PMDA approval: Heptavax-II (MSD K.K.) Bimmugen Bimmugen Injection 0.25 mL Bimmugen Injection 0.5 mL
Applicant: Kaketsuken [The Chemo-Sero-Therapeutic Research Institute]. Approval date: 2014-03-17. Type: Partial change. Active ingredient(s): Recombinant adsorbed hepatitis B vaccine (prepared from yeast).
Notes: Drugs with a revised dosage indicated for the prevention of perinatal hepatitis B virus infection (concomitant use with anti-Hepatitis B surface [HBs] human immunoglobulin). [Public knowledge-based application after PAFSC's preliminary assessment]